PMDA approval: (1) Skyrizi Intravenous Infusion 600 mg (2) Skyrizi for Subcutaneous Injection 360 mg Auto Dosers (3) Skyrizi for Subcutaneous Injection 180 mg Auto Dosers
Applicant: AbbVie GK. Approval date: 2024-06-24. Type: Partial change. Active ingredient(s): Risankizumab (genetical recombination).
Notes: 1) A drug with a new indication and a new dosage for the remission induction therapy of moderate to severe ulcerative colitis (for use only in patients who have not sufficiently responded to conventional treatments). (2) (3) Drugs with a new indication and a new dosage in an additional dosage form for the maintenance therapy for moderate to severe ulcerative colitis (for use only in patients who have not sufficiently responded to conventional treatments).